PMDA approval: Epclusa Combination Tablets
Applicant: Gilead Sciences, K.K. Approval date: 2022-08-24. Type: Partial change. Active ingredient(s): Sofosbuvir/ Velpatasvir.
Notes: A drug with a new indication and a new dosage for the improvement of viremia in patients with chronic hepatitis C or compensated cirrhosis type C who are naïve or have not previously been treated.